PMDA approval: Odefsey Combination Tablets
Applicant: Janssen Pharmaceutical K.K. Approval date: 2018-08-21. Type: New approval. Active ingredient(s): Rilpivirine hydrochloride /Emtricitabine/Tenofovir alafenamide fumarate.
Notes: A new combination drug with a new active ingredient indicated for the treatment of HIV-1 infection. [Orphan drug]